PMDA approval: Penles Tape 18 mg
Applicant: Nitto Denko Corporation. Approval date: 2013-06-14. Type: Partial change. Active ingredient(s): Lidocaine.
Notes: A drug with a new additional indication and a new dosage for the relief of pain during skin laser radiation therapy.